PMDA approval: Fintepla Oral Solution 2.2 mg/mL
Applicant: UCB Japan Co., Ltd. Approval date: 2022-09-26. Type: New approval. Active ingredient(s): Fenfluramine hydrochloride.
Notes: A drug with a new active ingredient indicated for use as an adjunctive therapy with other antiepileptic drugs to treat epileptic seizures in patients with Dravet syndrome who have not responded sufficiently to other antiepileptic drugs. [Orphan drug]